PMDA approval: Hemlibra s.c. 30 mg Hemlibra s.c. 60 mg Hemlibra s.c. 90 mg Hemlibra s.c. 105 mg Hemlibra s.c. 150 mg
Applicant: Chugai Pharmaceutical Co., Ltd. Approval date: 2018-12-21. Type: Partial change. Active ingredient(s): Emicizumab (genetical recombination).
Notes: Drugs with a new additional indication and a new dosage for the control of bleeding tendency in patients with blood coagulation factor VIII deficiency without blood coagulation factor VIII inhibitors.